PMDA approval: Xalkori Capsules 200 mg Xalkori Capsules 250 mg
Applicant: Pfizer Japan Inc. Approval date: 2017-05-18. Type: Partial change. Active ingredient(s): Crizotinib.
Notes: Drugs with a new additional indication for the treatment of unresectable advanced/relapsed ROS1 fusion gene-positive non-small-cell lung cancer. [Orphan drug]